PMDA approval: Linzess Tablets 0.25 mg
Applicant: Astellas Pharma Inc. Approval date: 2018-08-21. Type: Partial change. Active ingredient(s): Linaclotide.
Notes: A drug with a new additional indication for the treatment of chronic constipation (excluding constipation due to organic diseases).